PMDA approval: Januvia Tablets 25 mg Januvia Tablets 50 mg Januvia Tablets 100 mg (MSD K.K.) Glactiv Tablets 25 mg Glactiv Tablets 50 mg Glactiv Tablets 100 mg
Applicant: Ono Pharmaceutical Co., Ltd. Approval date: 2011-05-20. Type: Partial change. Active ingredient(s): Sitagliptin phosphate hydrate.
Notes: Drugs with a new additional indication for the treatment of type 2 diabetes mellitus in patients who have not responded sufficiently to alpha-glucosidase inhibitors along with diet and exercise therapies.